PMDA approval: Mitchga Vials 30 mg
Applicant: Maruho Co., Ltd. Approval date: 2024-03-26. Type: New approval. Active ingredient(s): Nemolizumab (genetical recombination).
Notes: A drug with a new indication and a new dosage for the treatment of prurigo nodularis in patients who have not responded sufficiently to conventional treatments, and a new additional pediatric dosage in an additional dosage form, indicated for the treatment of pruritus associated with atopic dermatitis in patients who have not responded sufficiently to conventional treatments.